PMDA approval: Prednisolone Tablets 5 mg "Takeda" Prednisolone Powder 1% "Takeda" (Takeda Pharmaceutical Company Limited) Predonine Tablets 5 mg
Applicant: Shionogi & Co., Ltd. Approval date: 2015-06-26. Type: Partial change. Active ingredient(s): Prednisolone.
Notes: Drugs with revised indications and a new dosage for the treatment of malignant lymphoma and similar diseases (closely-related diseases). [Expedited review]